PMDA approval: Ofev Capsules 100 mg Ofev Capsules 150 mg
Applicant: Nippon Boehringer Ingelheim Co., Ltd. Approval date: 2015-07-03. Type: New approval. Active ingredient(s): Nintedanib ethanesulfonate.
Notes: Drugs with a new active ingredient indicated for the treatment of idiopathic pulmonary fibrosis. [Orphan drug]